PMDA approval: Influenza HA Vaccine "Kitasatodaiichisankyo" Influenza HA Vaccine "S Hokken" Influenza HA Vaccine "Kitasatodaiichisankyo" Syringe
Applicant: Kitasato Daiichi Sankyo Vaccine Co., Ltd. Approval date: 2011-08-08. Type: Partial change. Active ingredient(s): Influenza HA Vaccine.
Notes: Drugs with a new pediatric dosage indicated for the prevention of influenza. [Expedited review]